PMDA approval: (1) Tocilizumab BS for Intravenous Infusion 80 mg “CT” (2) Tocilizumab BS for Intravenous Infusion 200 mg “CT” (3) Tocilizumab BS for Intravenous Infusion 400 mg “CT” (4) Tocilizumab BS 162 mg Syringes for S.C. Injection “CT” (5) Tocilizumab BS 162 mg Auto-Iinjector for S.C. Injection “CT”
Applicant: Celltrion Healthcare Japan K.K. Approval date: 2025-09-19. Type: New approval. Active ingredient(s): Tocilizumab (genetical recombination) [tocilizumab biosimilar 1].
Notes: (1) (2) (3) Biosimilars indicated for the following diseases: ・The treatment of rheumatoid arthritis (including the prevention of structural joint damage), polyarticular-course juvenile idiopathic arthritis, and systemic juvenile idiopathic arthritis, in patients who have not sufficiently responded to conventional treatments. ・The improvement of various symptoms and laboratory findings associated with Castleman's disease for whom resection of lymph nodes is not indicated. ・The treatment of cytokine release syndrome induced by the treatment of malignant tumor. (4) (5) Biosimilars indicated for the following disease ： ・The treatment of rheumatoid arthritis (including the prevention of structural joint damage) in patients who have not sufficiently responded to conventional treatments.